PMDA approval: Tracleer Tablets 62.5 mg
Applicant: Actelion Pharmaceuticals Japan Ltd. Approval date: 2015-08-24. Type: Partial change. Active ingredient(s): Bosentan hydrate.
Notes: A drug with a new additional indication for inhibiting development of digital ulcer in patients with systemic scleroderma (only for patients who currently have digital ulcers or have a history of digital ulcer.) [Orphan drug]